PMDA approval: Afinitor Tablets 5 mg
Applicant: Novartis Pharma K.K. Approval date: 2011-12-22. Type: Partial change. Active ingredient(s): Everolimus.
Notes: A drug with a new additional indication and a new dosage for the treatment of pancreatic neuroendocrine tumor.